PMDA approval: Vabysmo Solution for Intravitreal Injection 120 mg/mL
Applicant: Chugai Pharmaceutical Co., Ltd. Approval date: 2022-03-28. Type: New approval. Active ingredient(s): Faricimab (genetical recombination).
Notes: A drug with a new active ingredient indicated for the treatment of age-related macular degeneration associated with subfoveal choroidal neovascularization and diabetic macular edema.